PMDA approval: Beselna Cream 5%
Applicant: Mochida Pharmaceutical Co., Ltd. Approval date: 2011-11-25. Type: Partial change. Active ingredient(s): Imiquimod.
Notes: A drug with a new additional indication and a new dosage for the treatment of actinic keratosis (limited to the face or baldness).